PMDA approval: Golimumab BS 50 mg Syringe for S.C. Injection “F”
Applicant: Fuji Pharma Co., Ltd. Approval date: 2025-09-19. Type: New approval. Active ingredient(s): Golimumab (genetical recombination) [golimumab biosimilar 1].
Notes: A biosimilar indicated for the treatment of rheumatoid arthritis (including the prevention of structural joint damage) in patients who have not sufficiently responded to conventional treatments.